PMDA approval: [1] Sandostatin for S.C. Injection 50 μg Sandostatin for S.C. Injection 100 μg [2] Octreotide Acetate for S.C. Injection 50 μg “SANDOZ” Octreotide Acetate for S.C. Injection 100 μg “SANDOZ”
Applicant: [1] Novartis Pharma K.K. [2] Sandoz K.K. Approval date: 2020-08-21. Type: Partial change. Active ingredient(s): Octreotide acetate.
Notes: Drugs with a new additional indication and a new dosage for the treatment of hypoglycemia associated with congenital hyperinsulinism (for use only in patients who have not sufficiently responded to other pharmaceutical therapies ). [Public knowledge-based application after preliminary assessment by the Pharmaceutical Affairs and Food Sanitation Council (PAFSC)]